PMDA approval: Darzalex Intravenous Infusion 100 mg Darzalex Intravenous Infusion 400 mg
Applicant: Janssen Pharmaceutical K.K. Approval date: 2019-12-20. Type: Partial change. Active ingredient(s): Daratumumab (genetical recombination).
Notes: Drugs with a new dosage indicated for the treatment of multiple myeloma. [Orphan drug]